PMDA approval: Somatuline 120 mg for s.c. Injection
Applicant: Teijin Pharma Limited. Approval date: 2017-07-03. Type: Partial change. Active ingredient(s): Lanreotide acetate.
Notes: A drug with a new additional indication and a new dosage indicated for the treatment of neuroendocrine tumors of the pancreas and gastrointestinal tract.